PMDA approval: Simponi Subcutaneous Injection 50 mg Syringe
Applicant: Janssen Pharmaceutical K.K. Approval date: 2011-07-01. Type: New approval. Active ingredient(s): Golimumab (genetical recombination).
Notes: A drug with a new active ingredient indicated for the treatment of rheumatoid arthritis (including prevention of structural joint damage) in patients who have not sufficiently responded to conventional treatments.